PMDA approval: Pemetrexed for I.V. Infusion 100 mg “NK” Pemetrexed for I.V. Infusion 500 mg “NK” Pemetrexed for I.V. Infusion 800 mg “NK” Pemetrexed Solution for I.V. Infusion 100 mg “NK” Pemetrexed Solution for I.V. Infusion 500 mg “NK” Pemetrexed Solution for I.V. Infusion 800 mg “NK”
Applicant: Nippon Kayaku Co., Ltd. Approval date: 2023-03-27. Type: Partial change. Active ingredient(s): Pemetrexed sodium hemipentahydrate.
Notes: Drugs with a new indication and a new dosage for the preoperative adjuvant treatment for non-squamous non-small-cell lung cancer. [Expedited review]